PMDA approval: Ximency Combination Tablets
Applicant: Bristol-Myers Squibb K.K. Approval date: 2016-12-19. Type: New approval. Active ingredient(s): Daclatasvir hydrochloride/ Asunaprevir/ Beclabuvir hydrochloride.
Notes: A new combination drug with a new active ingredient indicated for the improvement of viremia in patients with chronic hepatitis C or compensated cirrhosis type C in serogroup 1 (genotype 1).